PMDA approval: (1) Comirnaty RTU Intramuscular Injection (2) Comirnaty RTU Intramuscular Injection for 1 person (3) Comirnaty Intramuscular Injection for 5 to 11 years old (4) Comirnaty Intramuscular Injection for 6 months to 4 years old
Applicant: Pfizer Japan Inc. Approval date: 2023-08-02. Type: Partial change. Active ingredient(s): (1) Tozinameran, Tozinameran and Famtozinameran, Tozinameran and Riltozinameran (2)-(4) Tozinameran, Tozinameran and Famtozinameran.
Notes: Drugs with a new indication, a new dosage, and other characteristics for the prevention of disease caused by SARS-CoV-2 infection (COVID-19). [Drug pertaining to Special Approval for Emergency]